PMDA approval: Spikevax Intramuscular Injection
Applicant: Moderna Japan Co., Ltd. Approval date: 2025-05-19. Type: Partial change. Active ingredient(s): mRNA encoding the spike protein of SARS-CoV-2.
Notes: A drug with a new additional pediatric dosage indicated for the prevention of disease caused by SARS-CoV-2 infection (COVID-19).